PMDA approval: Berinert S.C. Injection 2000
Applicant: CSL Behring K.K. Approval date: 2022-09-26. Type: New approval. Active ingredient(s): Human C1-inactivator.
Notes: A drug with a new route of administration indicated for the prophylaxis of acute attacks of hereditary angioedema. [Orphan drug]